PMDA approval: Utrogestan Vaginal Capsules 200 mg
Applicant: Fuji Pharma Co., Ltd. Approval date: 2016-01-22. Type: New approval. Active ingredient(s): Progesterone.
Notes: A drug with a new route of administration indicated for luteal support as part of assisted reproductive technology for infertile women.